PMDA approval: Dellegra Combination Tablets
Applicant: Sanofi K.K. Approval date: 2012-12-25. Type: New approval. Active ingredient(s): Fexofenadine hydrochloride/ pseudoephedrine hydrochloride.
Notes: A new combination drug indicated for the treatment of allergic rhinitis.